PMDA approval: Yervoy Injection 20 mg Yervoy Injection 50 mg
Applicant: Bristol-Myers Squibb K.K. Approval date: 2025-08-25. Type: Partial change. Active ingredient(s): Ipilimumab (genetical recombination).
Notes: Drugs with a new indication and new dosage for the treatment of unresectable, advanced or recurrent microsatellite instability-high (MSI-High) colorectal cancer. [Orphan drug]